PMDA approval: Pirespa Tablets 200 mg
Applicant: Shionogi & Co., Ltd. Approval date: 2008-10-16. Type: New approval. Active ingredient(s): Pirfenidone.
Notes: A drug containing a new active ingredient indicated for the treatment of idiopathic pulmonary fibrosis. [Orphan drug]